PMDA approval: Rituxan Injection 10 mg/mL
Applicant: Zenyaku Kogyo Co., Ltd. Approval date: 2015-05-26. Type: Partial change. Active ingredient(s): Rituximab (genetical recombination).
Notes: A drug with a new dosage for the treatment of CD20-positive, B-cell non-Hodgkin's lymphoma. [Orphan drug]